PMDA approval: (1) Bisono Tape 2 mg (2) Bisono Tape 4 mg (3) Bisono Tape 8 mg
Applicant: Toa Eiyo Ltd. Approval date: 2019-01-08. Type: New approval. Active ingredient(s): Bisoprolol.
Notes: (1) A drug with a new indication and a new dosage in an additional dosage form indicated for the treatment of tachycardiac atrial fibrillation. (2)(3) Drugs with a new additional indication, a new dosage, and other characteristics indicated for the treatment of tachycardiac atrial fibrillation.